PMDA approval: HyQvia 10％ S.C.Injection Set 5g/50mL HyQvia 10％ S.C.Injection Set 10g/100mL HyQvia 10％ S.C.Injection Set 20g/200mL
Applicant: Takeda Pharmaceutical Company Limited. Approval date: 2025-06-24. Type: Partial change. Active ingredient(s): pH4-treated acidic normal human immunoglobulin (subcutaneous injection) and Vorhyaluronidase alfa (genetical recombination).
Notes: Drugs with a new indication and a new dosage for inhibiting progression of motor disability due to chronic inflammatory demyelinating polyneuropathy and multifocal motor neuropathy (in the cases where patients show an improvement in their acute phase treatment).